PMDA approval: Botox Injection 50 Botox Injection 100
Applicant: GlaxoSmithKline K.K. Approval date: 2009-02-23. Type: Partial change. Active ingredient(s): Botulinum toxin type A.
Notes: Drugs with a new additional indication and a new dosage for the treatment of equinus deformity associated with lower limb spasticities in juvenile cerebral palsy in patients 2 years or older.